PMDA approval: Sotyktu Tablets 6 mg
Applicant: Bristol-Myers Squibb K.K. Approval date: 2022-09-26. Type: New approval. Active ingredient(s): Deucravacitinib.
Notes: A drug with a new active ingredient indicated for the treatment of plaque psoriasis, pustular psoriasis, and erythrodermic psoriasis in patients who have not responded sufficiently to conventional therapies.